PMDA approval: Prazaxa Capsules 75 mg Prazaxa Capsules 110 mg
Applicant: Nippon Boehringer Ingelheim Co., Ltd. Approval date: 2011-01-21. Type: New approval. Active ingredient(s): Dabigatran etexilate methanesulfonate.
Notes: Drugs with a new active ingredient indicated for prevention of ischemic stroke and systemic embolism in patients with nonvalvular atrial fibrillation.